PMDA approval: Vaxneuvance Aqueous Suspension Syringes
Applicant: MSD K.K. Approval date: 2022-09-26. Type: New approval. Active ingredient(s): Pneumococcal polysaccharides of serotypes 1, 3, 4, 5, 6A, 6B, 7F, 9V, 14, 18C, 19A, 19F, 22F, 23F, and 33F conjugated to CRM197 carrier protein.
Notes: A drug with a new active ingredient indicated for the prophylaxis of pneumococcal disease (serotypes 1, 3, 4, 5, 6A, 6B, 7F, 9V, 14, 18C, 19A, 19F, 22F, 23F, and 33F) in the elderly or in adults who are considered to be at a high risk of pneumococcal disease.